PMDA approval: Isovorin Injection 25 mg Isovorin Injection 100 mg
Applicant: Pfizer Japan Inc. Approval date: 2013-12-20. Type: Partial change. Active ingredient(s): Levofolinate calcium.
Notes: Drugs with a new additional indication and a new dosage for the treatment of unresectable pancreatic cancer. [Priority review], [Expedited review]